PMDA approval: Radiogardase Capsule 500 mg
Applicant: Nihon Medi-Physics Co., Ltd. Approval date: 2010-10-27. Type: New approval. Active ingredient(s): Iron(III)hexacyanoferrate(I I).
Notes: A drug with a new active ingredient indicated for the reduction of internal contamination with radioactive cesium. [Expedited review]